PMDA approval: Ustekinumab BS 45 mg Syringes for S.C. Injection “CT”
Applicant: Celltrion Healthcare Japan K.K. Approval date: 2025-03-27. Type: New approval. Active ingredient(s): Ustekinumab (genetical recombination) [ustekinumab biosimilar 3].
Notes: A biosimilar indicated for the treatment of the following disease in patients who have not sufficiently responded to conventional therapies: plaque psoriasis and psoriatic arthritis.